PMDA approval: Arikayce inhalation solution 590 mg
Applicant: Insmed GK. Approval date: 2021-03-23. Type: New approval. Active ingredient(s): Amikacin sulfate.
Notes: A drug with a new route of administration indicated for the treatment of nontuberculous mycobacterial pulmonary disease caused by amikacin-sensitive Mycobacterium avium complex (MAC).